PMDA approval: Tagrisso Tablets 40 mg Tagrisso Tablets 80 mg
Applicant: AstraZeneca K.K. Approval date: 2022-08-24. Type: Partial change. Active ingredient(s): Osimertinib mesilate.
Notes: Drugs with a new indication, a new dosage, and other characteristics for postoperative adjuvant treatment for epidermal growth factor receptor (EGFR) mutation-positive non-small cell lung cancer.